PMDA approval: (1) Stelara Intravenous Infusion 130 mg (2) Stelara Subcutaneous Injection 45 mg Syringe
Applicant: Janssen Pharmaceutical K.K. Approval date: 2017-03-30. Type: New approval. Active ingredient(s): Ustekinumab (genetical recombination).
Notes: (1) A drug with a new route of administration indicated for the induction therapy for moderate to severe active Crohn's disease (for use only in patients who have not sufficiently responded to conventional treatments). (2) A drug with a new indication and a new dosage for the maintenance therapy for moderate to severe active Crohn's disease (for use only in patients who have not sufficiently responded to conventional treatments).